PMDA approval: Istodax Injection 10 mg
Applicant: Celgene K.K. Approval date: 2017-07-03. Type: New approval. Active ingredient(s): Romidepsin.
Notes: A drug with a new active ingredient indicated for the treatment of relapsed or refractory peripheral T-cell lymphoma. [Orphan drug]